PMDA approval: G-Lasta Subcutaneous Injection 3.6 mg
Applicant: Kyowa Kirin Co., Ltd. Approval date: 2022-02-25. Type: Partial change. Active ingredient(s): Pegfilgrastim (genetical recombination).
Notes: A drug with a new indication and a new dosage for the mobilization of hematopoietic stem cell to the peripheral blood for allogenic peripheral blood stem cell transplantation.